PMDA approval: Januvia Tablets 25 mg Januvia Tablets 50 mg Januvia Tablets 100 mg (Banyu Pharmaceutical Co., Ltd.) Glactiv Tablets 25 mg Glactiv Tablets 50 mg Glactiv Tablets 100 mg
Applicant: Ono Pharmaceutical Co., Ltd. Approval date: 2009-10-16. Type: New approval. Active ingredient(s): Sitagliptin phosphate hydrate.
Notes: Drugs with a new active ingredient indicated for the treatment of type 2 diabetes mellitus (for use only in patients who do not sufficiently respond to any one of the following treatments): 1. Dietary therapy and/or exercise therapy only 2. Use of sulfonylureas in addition to dietary therapy and/or exercise therapy 3. Use of thiazolidinediones in addition to dietary therapy and/or exercise therapy 4. Use of biguanides in addition to dietary therapy and/or exercise therapy